PMDA approval: Skyrizi 150 mg for S.C. Injection Syringe 1 mL Skyrizi 150 mg for S.C. Injection Pen 1 mL Skyrizi 75 mg for S.C. Injection Syringe 0.83 mL
Applicant: AbbVie GK. Approval date: 2023-05-25. Type: Partial change. Active ingredient(s): Risankizumab (genetical recombination).
Notes: Drugs with a new indication and a new dosage for the treatment of palmoplantar pustulosis in patients who have not responded sufficiently to conventional therapies.